PMDA approval: Humira 40 mg for S.C. Injection Syringe 0.8 mL
Applicant: Abbott Japan Co., Ltd. Approval date: 2010-10-27. Type: Partial change. Active ingredient(s): Adalimumab (genetical recombination).
Notes: A drug with a new additional indication of ankylosing spondylitis (for use only in patients who have not sufficiently responded to conventional treatment). [Priority review]